PMDA approval: Randa Injection 10 mg/20 mL Randa Injection 25 mg/50 mL Randa Injection 50 mg/100 mL
Applicant: Nippon Kayaku Co., Ltd. Approval date: 2024-11-22. Type: Partial change. Active ingredient(s): Cisplatin.
Notes: Drugs with a new dosage indicated for the treatment of cervical cancer. [Expedited review]